PMDA approval: Cimzia 200 mg Syringe for S.C. Injection Cimzia 200 mg AutoClicks for S.C. Injection
Applicant: UCB Japan Co., Ltd. Approval date: 2019-12-20. Type: Partial change. Active ingredient(s): Certolizumab pegol (genetical recombination).
Notes: Drugs with new additional indications and a new dosage for the treatment of plaque psoriasis, psoriatic arthritis, pustular psoriasis, and erythrodermic psoriasis in patients who have not responded sufficiently to conventional therapies.